PMDA approval: Regnite Tablets 300 mg
Applicant: Astellas Pharma Inc. Approval date: 2012-01-18. Type: New approval. Active ingredient(s): Gabapentin enacarbil.
Notes: A drug with a new active ingredient indicated for the treatment of moderate to severe idiopathic restless legs syndrome.